PMDA approval: Kiklin Capsules 250 mg
Applicant: Astellas Pharma Inc. Approval date: 2016-02-29. Type: Partial change. Active ingredient(s): Bixalomer.
Notes: A drug with a new expanded indication for the improvement of hyperphosphatemia in patients with chronic kidney disease.